PMDA approval: Thymoglobuline for Intravenous Infusion 25 mg
Applicant: Sanofi-Aventis K.K. Approval date: 2008-07-16. Type: New approval. Active ingredient(s): Anti-human thymocyte immunoglobulin, Rabbit.
Notes: A drug containing a new active ingredient indicated for the treatment of moderate to very severe aplastic anemia, for use as pretreatment prior to hematopoietic stem cell transplantation, and for the treatment of acute graft-versus-host disease after hematopoietic stem cell transplantation. [Orphan drug]